PMDA approval: Xofluza Granules 2%
Applicant: Shionogi & Co., Ltd. Approval date: 2025-09-19. Type: Partial change. Active ingredient(s): Baloxavir marboxil.
Notes: A drug with a new additional pediatric dosage indicated for the treatment of influenza A or B virus infection.